PMDA approval: Kyprolis for Intravenous Injection10 mg Kyprolis for Intravenous Injection 40 mg
Applicant: Ono Pharmaceutical Co., Ltd. Approval date: 2016-07-04. Type: New approval. Active ingredient(s): Carfilzomib.
Notes: Drugs with a new active ingredient indicated for the treatment of relapsed or refractory multiple myeloma. [Orphan drug]